PMDA approval: Blopress Tablets 2 Blopress Tablets 4 Blopress Tablets 8 Blopress Tablets 12 (Teva Takeda Pharma Ltd.) Candesartan Tablets 2 mg “Aska” Candesartan Tablets 4 mg “Aska” Candesartan Tablets 8 mg “Aska” Candesartan Tablets 12 mg “Aska”
Applicant: Aska Pharmaceutical Co., Ltd. Approval date: 2019-05-22. Type: Partial change. Active ingredient(s): Candesartan cilexetil.
Notes: Drugs with a new additional pediatric dosage indicated for the treatment of hypertension. [Public knowledge-based application after preliminary assessment by the Pharmaceutical Affairs and Food Sanitation Council (PAFSC)]